PMDA approval: Harvoni Combination Tablets
Applicant: Gilead Sciences, Inc. Approval date: 2018-02-16. Type: Partial change. Active ingredient(s): Ledipasvir acetonate/Sofosbuvir.
Notes: A drug with a new additional indication for the improvement of viremia in patients with chronic hepatitis C or compensated cirrhosis type C in serogroup 2 (genotype 2).